PMDA approval: Naglazyme 5 mg for Intravenous Infusion
Applicant: AnGes MG, Inc. Approval date: 2008-03-28. Type: New approval. Active ingredient(s): Galsulfase (genetical recombination).
Notes: A drug containing a new active ingredient indicated for the treatment of mucopolysaccharidosis VI. [Orphan drug]